PMDA approval: Velexbru Tablets 80 mg
Applicant: Ono Pharmaceutical Co., Ltd. Approval date: 2020-08-21. Type: Partial change. Active ingredient(s): Tirabrutinib hydrochloride.
Notes: A drug with new indications for the treatment of Waldenström's macroglobulinemia and lymphoplasmacytic lymphoma. [Orphan drug]